PMDA approval: Aimovig Subcutaneous Injection 70 mg Pens
Applicant: Amgen K.K. Approval date: 2021-06-23. Type: New approval. Active ingredient(s): Erenumab (genetical recombination).
Notes: A drug with a new active ingredient indicated for the prevention of migraine attacks.